PMDA approval: Techne Phytate Kit
Applicant: PDRadiopharma Inc. Approval date: 2023-03-27. Type: Partial change. Active ingredient(s): Sodium phytate.
Notes: A drug with a new route of administration indicated for the sentinel lymph node mapping and lymphoscintigraphy in cervical cancer, uterine neoplasms, and head and neck cancer. A drug with a new indication for the sentinel lymph node mapping and lymphoscintigraphy in vulvar cancer. [Public knowledge-based application]